PMDA approval: Kostaive Intramuscular Injection
Applicant: Meiji Seika Pharma Co., Ltd. Approval date: 2024-09-13. Type: Partial change. Active ingredient(s): mRNA encoding the spike protein of SARS-CoV-2 (Omicron JN.1 variant).
Notes: A drug with a new indication, a new dosage, and other characteristics for the prevention of disease caused by SARS-CoV-2 infection (COVID-19). [Expedited review]